PMDA approval: Cystadane
Applicant: ReqMed Company, Ltd. Approval date: 2014-01-17. Type: New approval. Active ingredient(s): Betaine.
Notes: A drug with a new active ingredient indicated for the treatment of homocystinuria. [Orphan drug]